PMDA approval: Nevanac Ophthalmic Suspension 0.1%
Applicant: Alcon Japan Ltd. Approval date: 2010-10-27. Type: New approval. Active ingredient(s): Nepafenac.
Notes: A drug with a new active ingredient indicated for the treatment of postoperative inflammation in intraocular surgery.